PMDA approval: Kadcyla for Intravenous Infusion 100 mg Kadcyla for Intravenous Infusion 160 mg
Applicant: Chugai Pharmaceutical Co., Ltd. Approval date: 2020-08-21. Type: Partial change. Active ingredient(s): Trastuzumab emtansine (genetical recombination).
Notes: Drugs with a new indication and a new dosage for the postoperative adjuvant treatment for HER2-positive breast cancer.